PMDA approval: Toviaz Tablets 4 mg Toviaz Tablets 8 mg
Applicant: Pfizer Japan Inc. Approval date: 2022-09-26. Type: Partial change. Active ingredient(s): Fesoterodine fumarate.
Notes: Drugs with a new indication and a new additional pediatric dosage for the control of urination in patients with neurogenic bladder.